PMDA approval: Norditropin FlexPro Injection 5 mg Norditropin FlexPro Injection 10 mg Norditropin FlexPro Injection 15 mg Norditropin S Injection 10 mg
Applicant: Novo Nordisk Pharma Ltd. Approval date: 2017-11-30. Type: Partial change. Active ingredient(s): Somatropin (genetical recombination).
Notes: Drugs with a new additional indication for the treatment of short stature associated with Noonan syndrome with no epiphyseal closure.